PMDA approval: Adempas Tablets 0.5 mg Adempas Tablets 1.0 mg Adempas Tablets 2.5 mg
Applicant: Bayer Yakuhin, Ltd. Approval date: 2014-01-17. Type: New approval. Active ingredient(s): Riociguat.
Notes: Drugs with a new active ingredient indicated for the treatment of persistent/recurrent chronic thromboembolic pulmonary hypertension (CTEPH) after surgical treatment or inoperable CTEPH. [Orphan drug]